PMDA approval: Samsca Tablets 15 mg
Applicant: Otsuka Pharmaceutical Co., Ltd. Approval date: 2010-10-27. Type: New approval. Active ingredient(s): Tolvaptan.
Notes: A drug with a new active ingredient indicated for the treatment of fluid retention in heart failure patients who have not responded sufficiently to other diuretics to such as loop diuretics.